PMDA approval: Evoltra Intravenous Drip Infusion 20 mg
Applicant: Genzyme Japan K.K. Approval date: 2013-03-25. Type: New approval. Active ingredient(s): Clofarabine.
Notes: A drug with a new active ingredient for the treatment of recurrent or refractory acute lymphoblastic leukemia. [Orphan drug]